PMDA approval: Noxafil Tablets 100 mg Noxafil for Intravenous Infusion 300 mg
Applicant: MSD K.K. Approval date: 2020-01-23. Type: New approval. Active ingredient(s): Posaconazole.
Notes: Drugs with a new active ingredient indicated for the •prophylaxis of deep mycosis in hematopoietic stem cell transplantation recipients or patients with hematologic malignancy who are predicated to decrease neutrophil. •treatment of the following mycoses: Fusariosis, mucormycosis, coccidioidomycosis, chromoblastomycosis, and mycetoma.